PMDA approval: Braftovi Capsules 50 mg Braftovi Capsules 75 mg
Applicant: Ono Pharmaceutical Co., Ltd. Approval date: 2020-11-27. Type: Partial change. Active ingredient(s): Encorafenib.
Notes: Drugs with a new indication and a new dosage for the treatment of unresectable, advanced or recurrent BRAF mutation-positive colorectal cancer that has progressed after cancer chemotherapy. [Priority review]